PMDA approval: Aselend Injection 100 μg
Applicant: Fujimoto Pharmaceutical Corporation. Approval date: 2019-03-26. Type: New approval. Active ingredient(s): Sodium selenite.
Notes: A drug with a new active ingredient indicated for the treatment of hyposelenemia.